PMDA approval: Solu-Cortef Injection 100 mg Solu-Cortef for Intravenous Use 250 mg Solu-Cortef for Intravenous Use 500 mg
Applicant: Pfizer Japan Inc. Approval date: 2015-05-26. Type: Partial change. Active ingredient(s): Hydrocortisone sodium succinate.
Notes: Drugs with a new additional indication and a new dosage for the treatment of bronchial asthma. [Public knowledge-based application after PAFSC's preliminary assessment]